PMDA approval: Imidafenacin Uritos Tablets 0.1 mg (Kyorin Pharmaceutical Co., Ltd.) Staybla Tablets 0.1 mg
Applicant: Ono Pharmaceutical Co., Ltd. Approval date: 2007-04-18. Type: New approval. Active ingredient(s): Imidafenacin.
Notes: Drugs containing a new active ingredient indicated for the treatment of micturition urgency, pollakiuria, and urge incontinence associated with overactive bladder.